PMDA approval: Gonalef for Subcutaneous Injection 150
Applicant: Merck Serono Co., Ltd. Approval date: 2011-05-20. Type: Partial change. Active ingredient(s): Follitropin alfa (genetical recombination).
Notes: A drug with a new additional indication and a new dosage for induction of ovulation in patients with anovulation and infrequent ovulation associated with hypothalamic-pituitary dysfunction or polycystic ovarian syndrome.